PMDA approval: Daklinza Tablets 60 mg (Bristol-Myers K.K.) Sunvepra Capsules 100 mg
Applicant: Bristol-Myers K.K. Approval date: 2014-07-04. Type: New approval. Active ingredient(s): Daclatasvir hydrochloride Asunaprevir.
Notes: Drugs with new active ingredients indicated for the improvement of viremia in patients with chronic hepatitis C or compensated cirrhosis type C in serogroup 1 (genotype 1) who are untreated and ineligible for, or are intolerant to therapy including interferon; or who did not respond to therapy including interferon. [Priority review]